PMDA approval: Tamiflu Dry Syrup 3%
Applicant: Chugai Pharmaceutical Co., Ltd. Approval date: 2017-03-24. Type: Partial change. Active ingredient(s): Oseltamivir phosphate.
Notes: A drug with a new additional dosage for newborns and infants for the treatment of influenza A or B virus infection. [Public knowledge-based application after PAFSC's preliminary assessment]